PMDA approval: Inderal Tablets 10 mg Inderal Tablets 20 mg
Applicant: AstraZeneca K.K. Approval date: 2012-05-25. Type: Partial change. Active ingredient(s): Propranolol hydrochloride.
Notes: Drugs with new additional pediatric dosages for extrasystole (supraventricular/ventricular), the prevention of paroxysmal tachycardia, atrial fibrillation with rapid ventricular response (bradycardiac effect), sinus tachycardia, atrial fibrillation, and the prevention of paroxysmal atrial fibrillation. [Public knowledge-based application after PAFSC's preliminary assessment]